PMDA approval: Emla Cream
Applicant: Sato Pharmaceutical Co., Ltd. Approval date: 2012-01-18. Type: New approval. Active ingredient(s): Lidocaine/propitocaine.
Notes: A new combination drug with a new active ingredient indicated for the relief of pain in skin laser radiation therapy.